PMDA approval: Vyvanse Capsules 20 mg Vyvanse Capsules 30 mg
Applicant: Shionogi & Co., Ltd. Approval date: 2019-03-26. Type: New approval. Active ingredient(s): Lisdexamfetamine mesilate.
Notes: Drugs with a new active ingredient indicated for the treatment of pediatric attention deficit/hyperactivity disorder (AD/HD).